PMDA approval: Darbepoetin Alfa BS 5 μg Syringe for Injection “Sanwa” Darbepoetin Alfa BS 10 μg Syringe for Injection “Sanwa” Darbepoetin Alfa BS 15 μg Syringe for Injection “Sanwa” Darbepoetin Alfa BS 20 μg Syringe for Injection “Sanwa” Darbepoetin Alfa BS 30 μg Syringe for Injection “Sanwa” Darbepoetin Alfa BS 40 μg Syringe for Injection “Sanwa” Darbepoetin Alfa BS 60 μg Syringe for Injection “Sanwa” Darbepoetin Alfa BS 120 μg Syringe for Injection “Sanwa” Darbepoetin Alfa BS 180 μg Syringe for Injection “Sanwa”
Applicant: Sanwa Kagaku Kenkyusho Co., Ltd. Approval date: 2019-09-20. Type: New approval. Active ingredient(s): Darbepoetin alfa (genetical recombination) [darbepoetin alfa biosimilar 2].
Notes: Follow-on biologics indicated for the treatment of nephrogenic anaemia.